PMDA approval: Enaroy Tablets 2 mg Enaroy Tablets 4 mg
Applicant: Japan Tobacco Inc. Approval date: 2020-09-25. Type: New approval. Active ingredient(s): Enarodustat.
Notes: Drugs with a new active ingredient indicated for the treatment of renal anemia.